PMDA approval: Actemra 80 mg for Intravenous Infusion Actemra 200 mg for Intravenous Infusion Actemra 400 mg for Intravenous Infusion
Applicant: Chugai Pharmaceutical Co., Ltd. Approval date: 2019-03-26. Type: Partial change. Active ingredient(s): Tocilizumab (genetical recombination).
Notes: Drugs with a new additional indication and a new dosage for the treatment of cytokine release syndrome induced by tumor-specific T-cell infusion therapy. [Expedited review]